PMDA approval: Onoact 50 for injection
Applicant: Ono Pharmaceutical Co., Ltd. Approval date: 2013-11-22. Type: Partial change. Active ingredient(s): Landiolol hydrochloride.
Notes: A drug with a new additional indication and a new dosage for the treatment of tachyarrhythmia including atrial fibrillation/flutter in patients with low cardiac function.